PMDA approval: Atedio Combination Tab.
Applicant: Ajinomoto Pharmaceutical Co., Ltd. Approval date: 2014-03-24. Type: New approval. Active ingredient(s): Valsartan/cilnidipine.
Notes: A new combination drug indicated for the treatment of hypertension.